PMDA approval: Rexulti Tablets 1 mg Rexulti Tablets 2 mg Rexulti OD Tablets 0.5 mg Rexulti OD Tablets 1 mg Rexulti OD Tablets 2 mg
Applicant: Otsuka Pharmaceutical Co., Ltd. Approval date: 2023-12-22. Type: Partial change. Active ingredient(s): Brexpiprazole.
Notes: Drugs with a new indication and a new dosage for the treatment of depression (for use only in patients who have not sufficiently responded to conventional treatments).